PMDA approval: Fludara I.V.Infusion 50 mg
Applicant: Sanofi K.K. Approval date: 2022-06-20. Type: Partial change. Active ingredient(s): Fludarabine phosphate.
Notes: A drug with a new indication and a new dosage for the treatment of relapsed or refractory acute myeloid leukemia. [Public knowledge-based application after PAFSC's preliminary assessment]